PMDA approval: Bevacizumab BS Intravenous Infusion 100 mg [Pfizer] Bevacizumab BS Intravenous Infusion 400 mg [Pfizer]
Applicant: Pfizer Japan Inc. Approval date: 2019-06-18. Type: New approval. Active ingredient(s): Bevacizumab (genetical recombination) [bevacizumab biosimilar 1].
Notes: Follow-on biologics indicated for the treatment of unresectable advanced or recurrent colorectal cancer.